PMDA approval: Opdivo Intravenous Infusion 20 mg Opdivo Intravenous Infusion 100 mg Opdivo Intravenous Infusion 120 mg Opdivo Intravenous Infusion 240 mg
Applicant: Ono Pharmaceutical Co., Ltd. Approval date: 2023-11-24. Type: Partial change. Active ingredient(s): Nivolumab (genetical recombination).
Notes: Drugs with a new indication and a new dosage for the treatment of malignant mesothelioma (excluding malignant pleural mesothelioma). [Orphan drug]